PMDA approval: Ticovac Suspension Liquid for Intramuscular Injection 0.5 mL Ticovac Junior Suspension Liquid for Intramuscular Injection 0.25 mL
Applicant: Pfizer Japan Inc. Approval date: 2024-03-26. Type: New approval. Active ingredient(s): Inactivated tick-borne encephalitis virus.
Notes: Drugs with a new active ingredient indicated for the prevention of tick-borne encephalitis.